PMDA approval: Norspan Tape 5 mg Norspan Tape 10 mg Norspan Tape 20 mg
Applicant: Mundipharma K.K. Approval date: 2011-02-23. Type: New approval. Active ingredient(s): Buprenorphine.
Notes: Drugs with a new active ingredient indicated for analgesia of chronic pain associated with osteoarthritis and lumbago which cannot be managed by treatments with non-opioid analgesics.